PMDA approval: Eylea Intravitreal Injection 40 mg/mL
Applicant: Bayer Yakuhin, Ltd. Approval date: 2022-09-26. Type: Partial change. Active ingredient(s): Aflibercept (genetical recombination).
Notes: A drug with a new indication and a new dosage for the treatment of retinopathy of prematurity.